PMDA approval: MabCampath I.V. Infusion 30 mg
Applicant: Sanofi K.K. Approval date: 2020-12-25. Type: Partial change. Active ingredient(s): Alemtuzumab (genetical recombination).
Notes: A drug with a new indication and a new dosage for a conditioning regimen prior to allogeneic hematopoietic stem cell transplantation. [Orphan drug]